PMDA approval: Ancaron Tablets 100
Applicant: Sanofi-Aventis K.K. Approval date: 2010-09-10. Type: Partial change. Active ingredient(s): Amiodarone hydrochloride.
Notes: A drug with a new additional indication for the treatment of arterial fibrillation with cardiac failure (impaired cardiac function).